PMDA approval: Poteligeo Injection 20 mg
Applicant: Kyowa Hakko Kirin Co., Ltd. Approval date: 2014-12-18. Type: Partial change. Active ingredient(s): Mogamulizumab (genetical recombination).
Notes: A drug with a revised indication and a new dosage for the treatment of CCR4-positive adult T-cell leukemia/lymphoma. [Expedited review]